PMDA approval: Aimmugen
Applicant: Kaketsuken [The Chemo-Sero-Therapeutic Research Institute]. Approval date: 2013-03-15. Type: Partial change. Active ingredient(s): Freeze-dried inactivated tissue culture hepatitis A vaccine.
Notes: A drug with a new pediatric dosage. The drug is indicated for the prevention of hepatitis A.